PMDA approval: Samsca Tablets 7.5 mg Samsca Tablets 15 mg Samsca Tablets 30 mg Samsca OD Tablets 7.5 mg Samsca OD Tablets 15 mg Samsca OD Tablets 30 mg Samsca Granules 1%
Applicant: Otsuka Pharmaceutical Co., Ltd. Approval date: 2020-06-29. Type: Partial change. Active ingredient(s): Tolvaptan.
Notes: Drugs with a new indication and a new dosage for the improvement of hyponatraemia in patients with syndrome of inappropriate secretion of antidiuretic hormone (SIADH). [Orphan drug]